PMDA approval: Tramal Capsules 25 mg Tramal Capsules 50 mg
Applicant: Nippon Shinyaku Co., Ltd. Approval date: 2010-07-23. Type: New approval. Active ingredient(s): Tramadol hydrochloride.
Notes: Drugs with a new route of administration indicated for analgesia in various types of cancer with mild to moderate pain.